PMDA approval: Xalacom Combination Eye Drops
Applicant: Pfizer Japan Inc. Approval date: 2010-01-20. Type: New approval. Active ingredient(s): Latanoprost/timolol maleate.
Notes: A new combination drug indicated for the treatment of glaucoma and ocular hypertension.